PMDA approval: Spiriva 2.5 μg Respimat 60 puffs
Applicant: Nippon Boehringer Ingelheim Co., Ltd. Approval date: 2014-11-18. Type: Partial change. Active ingredient(s): Tiotropium bromide hydrate.
Notes: A drug with a new additional indication for the relief of symptoms secondary to airway obstructive disorders in bronchial asthma (for use only in patients with the severe persistent type).